PMDA approval: Stivarga tablets 40 mg
Applicant: Bayer Yakuhin, Ltd. Approval date: 2013-03-25. Type: New approval. Active ingredient(s): Regorafenib hydrate.
Notes: A drug with a new active ingredient indicated for the treatment of unresectable advanced or recurrent colorectal cancer. [Priority review]